PMDA approval: Lunabell Tablets LD
Applicant: Nobelpharma Co., Ltd. Approval date: 2022-03-11. Type: Partial change. Active ingredient(s): Norethisterone/ Ethinylestradiol.
Notes: A drug with a new indication and a new dosage for adjusting the start timing of controlled ovarian stimulation in assisted reproductive technology. [Public knowledge-based application]